PMDA approval: Tecentriq Intravenous Infusion 1200 mg
Applicant: Chugai Pharmaceutical Co., Ltd. Approval date: 2022-05-26. Type: Partial change. Active ingredient(s): Atezolizumab (genetical recombination).
Notes: A drug with a new indication and a new dosage for the postoperative adjuvant treatment for PD-L1-positive non-small cell lung cancer.